PMDA approval: Rinvoq Tablets 7.5 mg Rinvoq Tablets 15 mg
Applicant: AbbVie GK. Approval date: 2022-05-26. Type: Partial change. Active ingredient(s): Upadacitinib hydrate.
Notes: Drugs with a new indication and a new dosage for the treatment of ankylosing spondylitis in patients who have not responded sufficiently to conventional treatments.